PMDA approval: Bevacizumab BS for Intravenous Infusion 100 mg “CTNK” Bevacizumab BS for Intravenous Infusion 400 mg “CTNK”
Applicant: Nippon Kayaku Co., Ltd. Approval date: 2022-09-26. Type: New approval. Active ingredient(s): Bevacizumab (genetical recombination) [bevacizumab biosimilar 4].
Notes: Follow-on biologics indicated for the treatment of unresectable advanced or recurrent colon or rectal cancer, unresectable advanced or recurrent non-squamous non-small cell lung cancer, and unresectable or recurrent breast cancer.